PMDA approval: Gemzar Injection 200 mg Gemzar Injection 1 g
Applicant: Eli Lilly Japan K.K. Approval date: 2008-11-25. Type: Partial change. Active ingredient(s): Gemcitabine hydrochloride.
Notes: Drugs with a new additional indication for the treatment of urothelial carcinoma.